PMDA approval: Hernexeos Tablets 60 mg
Applicant: Nippon Boehringer Ingelheim Co., Ltd. Approval date: 2025-09-19. Type: New approval. Active ingredient(s): Zongertinib.
Notes: A drug with a new active ingredient indicated for the treatment of unresectable advanced or recurrent HER2 (ERBB2 ) mutation-positive non-small cell lung cancer that has progressed after cancer chemotherapy. [Orphan drug]